PMDA approval: BeneFIX Intravenous 250 BeneFIX Intravenous 500 BeneFIX Intravenous 1000 BeneFIX Intravenous 2000
Applicant: Wyeth K.K. Approval date: 2009-10-16. Type: New approval. Active ingredient(s): Nonacog alfa (genetical recombination).
Notes: Drugs with a new active ingredient indicated for inhibition of bleeding tendency in patients with hemophilia B (congenital blood coagulation factor IX deficiency). [Orphan drug]